PMDA approval: Keytruda Injection 20 mg Keytruda Injection 100 mg
Applicant: MSD K.K. Approval date: 2019-12-20. Type: Partial change. Active ingredient(s): Pembrolizumab (genetical recombination).
Notes: 1) Drugs with a new indication and a new dosage for the treatment of unresectable or metastatic renal cell carcinoma. 2) Drugs with a new indication for the treatment of recurrent or metastatic head and neck cancer.